PMDA approval: Ninlaro Capsules 2.3 mg Ninlaro Capsules 3 mg Ninlaro Capsules 4 mg
Applicant: Takeda Pharmaceutical Company Limited. Approval date: 2017-03-30. Type: New approval. Active ingredient(s): Ixazomib citrate.
Notes: Drugs with a new active ingredient indicated for the treatment of relapsed or refractory multiple myeloma. [Orphan drug]